PMDA approval: Vynmac Capsules 61 mg
Applicant: Pfizer Japan Inc. Approval date: 2021-09-27. Type: New approval. Active ingredient(s): Tafamidis.
Notes: A drug with a new active ingredient indicated for the treatment of transthyretin cardiac amyloidosis (wild-type and hereditary).